PMDA approval: Jakavi Tablets 5 mg Jakavi Tablets 10 mg Jakavi Oral Solution for Pediatric 0.5%
Applicant: Novartis Pharma K.K. Approval date: 2024-09-24. Type: Partial change. Active ingredient(s): Ruxolitinib phosphate.
Notes: Drugs with a new indication and a new additional pediatric dosage and a drug with a new indication and a new additional pediatric dosage in an additional dosage form for the treatment of graft-versus-host disease after hematopoietic stem cell transplantation (for use when steroid drugs are not sufficiently effective). [Orphan drug]